PMDA approval: E Keppra Tablets 250 mg E Keppra Tablets 500 mg
Applicant: UCB Japan Co., Ltd. Approval date: 2010-07-23. Type: New approval. Active ingredient(s): Levetiracetam.
Notes: Drugs with a new active ingredient indicated for use as an adjunctive therapy with other antiepileptic drugs to treat partial seizures (including secondary generalized seizures) in patients with epilepsy who have not responded sufficiently to other antiepileptic drugs.